PMDA approval: Xtandi Capsules 40 mg
Applicant: Astellas Pharma Inc. Approval date: 2014-03-24. Type: New approval. Active ingredient(s): Enzalutamide.
Notes: A drug with a new active ingredient indicated for the treatment of castration-resistant prostate cancer. [Priority review]